PMDA approval: Onon Drysyrup 10%
Applicant: Ono Pharmaceutical Co., Ltd. Approval date: 2011-12-22. Type: Partial change. Active ingredient(s): Pranlukast hydrate.
Notes: A drug with a new additional indication for the treatment of allergic rhinitis in pediatric patients.